PMDA approval: Brazaves Capsules 100 mg
Applicant: Actelion Pharmaceuticals Japan Ltd. Approval date: 2012-03-30. Type: New approval. Active ingredient(s): Miglustat.
Notes: A drug with a new active ingredient indicated for the treatment of Niemann-Pick disease Type C. [Orphan drug]